PMDA approval: Treakisym Injection 25 mg Treakisym Injection 100 mg
Applicant: SymBio Pharmaceuticals Limited. Approval date: 2021-03-23. Type: Partial change. Active ingredient(s): Bendamustine hydrochloride.
Notes: Drugs with a new indication and a new dosage for the treatment of relapsed or refractory diffuse large B-cell lymphoma. [Expedited review]